PMDA approval: Zoladex LA 10.8 mg depot
Applicant: AstraZeneca K.K. Approval date: 2014-03-17. Type: Partial change. Active ingredient(s): Goserelin acetate.
Notes: A drug with a new additional indication for the treatment of premenopausal breast cancer.